PMDA approval: Durotep MT Patch 2.1 mg Durotep MT Patch 4.2 mg Durotep MT Patch 8.4 mg Durotep MT Patch 12.6 mg Durotep MT Patch 16.8 mg
Applicant: Janssen Pharmaceutical K.K. Approval date: 2008-03-19. Type: New approval. Active ingredient(s): Fentanyl.
Notes: Drugs with a new dosage form indicated for pain relief in cancer patients with moderate to severe pain.